PMDA approval: [1] Abilify Tablets 3 mg Abilify Tablets 6 mg Abilify Tablets 12 mg Abilify Powder 1% Abilify Oral Solution 0.1% [2] Abilify OD Tablets 3 mg Abilify OD Tablets 6 mg Abilify OD Tablets 12 mg Abilify OD Tablets 24 mg
Applicant: Otsuka Pharmaceutical Co., Ltd. Approval date: 2012-01-18. Type: Partial change. Active ingredient(s): Aripiprazole.
Notes: Drugs with a new additional indication and a new dosage in new dosage forms for the improvement of manic symptoms in patients with bipolar disorder.